PMDA approval: P-Tol Chewable Tab. 250 mg P-Tol Chewable Tab. 500 mg
Applicant: Kissei Pharmaceutical Co., Ltd. Approval date: 2015-09-28. Type: New approval. Active ingredient(s): Sucroferric oxyhydroxide.
Notes: Drugs with a new active ingredient indicated for the improvement of hyperphosphatemia in patients with chronic kidney disease who are receiving dialysis.